PMDA approval: Xeomin 50 Units for Intramuscular Injection Xeomin 100 Units for Intramuscular Injection Xeomin 200 Units for Intramuscular Injection
Applicant: Teijin Pharma Limited. Approval date: 2020-06-29. Type: New approval. Active ingredient(s): Incobotulinumtoxin A.
Notes: Drugs with a new active ingredient indicated for the treatment of upper limb spasticity.